PMDA approval: INOflo for Inhalation 800 ppm
Applicant: INO therapeutics LLC. Approval date: 2008-07-16. Type: New approval. Active ingredient(s): Nitric oxide.
Notes: A drug containing a new active ingredient indicated for the treatment of hypoxic respiratory failure (HRF) with concurrent pulmonary hypertension in neonates. [Orphan drug]